PMDA approval: Olumiant Tablets 2 mg Olumiant Tablets 4 mg
Applicant: Eli Lilly Japan K.K. Approval date: 2022-06-20. Type: Partial change. Active ingredient(s): Baricitinib.
Notes: Drugs with a new indication and a new dosage for the treatment of alopecia areata (for use only in patients with intractable and extensive alopecia areata).